PMDA approval: Fycompa Tablets 2 mg Fycompa Tablets 4 mg
Applicant: Eisai Co., Ltd. Approval date: 2016-03-28. Type: New approval. Active ingredient(s): Perampanel hydrate.
Notes: Drugs with a new active ingredient indicated for use as an adjunctive therapy with other antiepileptic drugs to treat partial seizures (including secondary generalized seizures) and tonic-clonic seizures in patients with epilepsy who have not responded sufficiently to other antiepileptic drugs.